PMDA approval: Acoalan Injection 600
Applicant: Kyowa Hakko Kirin Co., Ltd. Approval date: 2015-07-03. Type: New approval. Active ingredient(s): Antithrombin gamma (genetical recombination).
Notes: A drug with a new active ingredient indicated for the treatment of thrombophilia due to congenital antithrombin (AT) deficiency (CAD) and disseminated intravascular coagulation (DIC) accompanied by a decrease in AT.